PMDA approval: Retevmo Capsules 40 mg Retevmo Capsules 80 mg
Applicant: Eli Lilly Japan K.K. Approval date: 2021-09-27. Type: New approval. Active ingredient(s): Selpercatinib.
Notes: Drugs with a new active ingredient indicated for the treatment of RET fusion gene-positive unresectable advanced or recurrent non-small cell lung cancer. [Orphan drug]